PMDA approval: Glubes Combination Tab.
Applicant: Kissei Pharmaceutical Co., Ltd. Approval date: 2011-04-22. Type: New approval. Active ingredient(s): Mitiglinide calcium hydrate/voglibose.
Notes: A new combination drug indicated for the treatment of type 2 diabetes mellitus (only when a concomitant use of mitiglinide calcium hydrate with voglibose is deemed appropriate).